PMDA approval: Lokelma 5 g Powder for Suspension (Single-dose package) Lokelma 10 g Powder for Suspension (Single-dose package)
Applicant: AstraZeneca K.K. Approval date: 2020-03-25. Type: New approval. Active ingredient(s): Sodium zirconium cyclosilicate hydrate.
Notes: Drugs with a new active ingredient indicated for the treatment of hyperkalaemia.